PMDA approval: Noxafil Tablets 100 mg Noxafil for Intravenous Infusion 300 mg
Applicant: MSD K.K. Approval date: 2021-09-27. Type: Partial change. Active ingredient(s): Posaconazole.
Notes: Drugs with a new indication for the treatment of invasive aspergillosis.